PMDA approval: Actemra 162 mg Syringe for SC Injection Actemra 162 mg Auto-Injector for SC Injection
Applicant: Chugai Pharmaceutical Co., Ltd. Approval date: 2013-03-25. Type: New approval. Active ingredient(s): Tocilizumab (genetical recombination).
Notes: Drugs with a new route of administration indicated for the treatment of rheumatoid arthritis (including prevention of structural joint damage) in patients who have not sufficiently responded to conventional treatments.